PMDA approval: Solu-Medrol for Intravenous Use 40 mg Solu-Medrol for Intravenous Use 125 mg Solu-Medrol for Intravenous Use 500 mg Solu-Medrol for Intravenous Use 1000 mg
Applicant: Pfizer Japan Inc. Approval date: 2023-09-25. Type: Partial change. Active ingredient(s): Methylprednisolone sodium succinate.
Notes: Drugs with a new indication and a new dosage for the treatment of the acute phase of Kawasaki's disease (for patients with severe Kawasaki's disease who have the risk of developing coronary artery disorder). [Public knowledge-based application after PAFSC's preliminary assessment]